PMDA approval: Gonalef for Subcutaneous Injection 75 Gonalef for Subcutaneous Injection 150 Gonalef for Subcutaneous Injection Pen 300 Gonalef for Subcutaneous Injection Pen 450 Gonalef for Subcutaneous Injection Pen 900
Applicant: Merck Serono Co., Ltd. Approval date: 2019-02-21. Type: Partial change. Active ingredient(s): Follitropin alfa (genetical recombination).
Notes: Drugs with changes in dosage regimen indicated for the induction of spermatogenesis in male hypogonadotropic hypogonadism (MHH) and with a new additional indication and a new dosage for controlled ovarian stimulation in assisted reproductive technology.